PMDA approval: Humira 40 mg for S.C. Injection Syringe 0.4 mL Humira 80 mg for S.C. Injection Syringe 0.8 mL Humira 40 mg for S.C. Injection in pre-filled pen 0.4 mL Humira 80 mg for S.C. Injection in pre-filled pen 0.8 mL
Applicant: AbbVie GK. Approval date: 2020-11-27. Type: Partial change. Active ingredient(s): Adalimumab (genetical recombination).
Notes: Drugs with a new indication and a new dosage for the treatment of pyoderma gangrenosum. [Orphan drug]